PMDA approval: Tecentriq Intravenous Infusion 1200 mg
Applicant: Chugai Pharmaceutical Co., Ltd. Approval date: 2020-12-25. Type: Partial change. Active ingredient(s): Atezolizumab (genetical recombination).
Notes: A drug with a new dosage indicated for the treatment of unresectable advanced or recurrent non-small cell lung cancer.